PMDA approval: Enefluid Injection
Applicant: Otsuka Pharmaceutical Factory, Inc. Approval date: 2020-09-25. Type: New approval. Active ingredient(s): N/A for this combination drug.
Notes: A combination prescription drug with similar formulations indicated for the supplementation of amino acids, electrolytes, calories, fatty acids, water-soluble vitamin, and water in the following conditions. •When patients with insufficient oral intake and mild hypoproteinemia or mild undernutrition. •When patients in the perioperative period